PMDA approval: Cresemba Capsules 100 mg Cresemba for I.V. Infusion 200 mg
Applicant: Asahi Kasei Pharma Corporation. Approval date: 2022-12-23. Type: New approval. Active ingredient(s): Isavuconazonium sulfate.
Notes: Drugs with a new active ingredient indicated for the treatment of the following mycoses: • Aspergillosis (invasive aspergillosis, chronic progressive pulmonary aspergillosis, simple pulmonary aspergilloma) • Mucormycosis • Cryptococcosis (pulmonary cryptococcosis, disseminated cryptococcosis [including cryptococcal meningitis])